PMDA approval: Lotriga Granular Capsule 2 g
Applicant: Takeda Pharmaceutical Company Limited. Approval date: 2012-09-28. Type: New approval. Active ingredient(s): omega-3-acid ethyl esters.
Notes: A drug with a new active ingredient indicated for the treatment of hyperlipidaemia.